PMDA approval: Quintovac Aqueous Suspension Injection
Applicant: KM Biologics Co., Ltd. Approval date: 2023-09-25. Type: New approval. Active ingredient(s): Protective antigen of Bordetella pertussis Diphtheria toxoid Tetanus toxoid Inactivated poliovirus types 1-3 (Sabin strain) Haemophilus influenzae type b polysaccharide-tetanus toxoid conjugate.
Notes: A new combination drug with a new active ingredient indicated for the prevention of pertussis, diphtheria, tetanus, acute poliomyelitis, and infections caused by Haemophilus influenzae type b.